PMDA approval: Lumakras Tablets 120 mg
Applicant: Amgen K.K. Approval date: 2025-09-19. Type: Partial change. Active ingredient(s): Sotorasib.
Notes: A drug with a new indication and a new dosage for the treatment of unresectable advanced or recurrent KRAS G12C mutation-positive colorectal cancer that has progressed after cancer chemotherapy.